PMDA approval: Flagyl Oral Tablet 250 mg Flagyl Vaginal Tablet 250 mg
Applicant: Shionogi & Co., Ltd. Approval date: 2012-03-21. Type: Partial change. Active ingredient(s): Metronidazole.
Notes: Drugs with a new indication and new dosages for the treatment of bacterial vaginosis. [Public knowledge-based application after PAFSC's preliminary assessment]